PMDA approval: Vizimpro Tablets 15 mg Vizimpro Tablets 45 mg
Applicant: Pfizer Japan Inc. Approval date: 2019-01-08. Type: New approval. Active ingredient(s): Dacomitinib hydrate.
Notes: Drugs with a new active ingredient indicated for the treatment of inoperable or recurrent non-small cell lung cancer with EGFR gene mutation. [Priority review]